PMDA approval: Glanatec Ophthalmic Solution 0.4%
Applicant: Kowa Company, Ltd. Approval date: 2014-09-26. Type: New approval. Active ingredient(s): Ripasudil hydrochloride hydrate.
Notes: A drug with a new active ingredient indicated for the treatment of glaucoma or ocular hypertension in patients who have not sufficiently responded to other therapeutic drugs for glaucoma or who are unable to use them.